PMDA approval: Romiplate for S.C. Injection 250 μg
Applicant: Kyowa Hakko Kirin Co., Ltd. Approval date: 2011-01-21. Type: New approval. Active ingredient(s): Romiplostim (genetical recombination).
Notes: A drug with a new active ingredient indicated for the treatment of chronic idiopathic thrombocytopenic purpura. [Orphan drug]